PMDA approval: Endoxan for Injection 100 mg Endoxan for Injection 500 mg Endoxan Tablets 50 mg
Applicant: Shionogi & Co., Ltd. Approval date: 2021-08-25. Type: Partial change. Active ingredient(s): Cyclophosphamide hydrate.
Notes: Drugs with a new indication and a new dosage for the treatment of systemic AL amyloidosis. [Expedited review]